PMDA approval: Bosulif Tablets 100 mg
Applicant: Pfizer Japan Inc. Approval date: 2020-06-29. Type: Partial change. Active ingredient(s): Bosutinib hydrate.
Notes: A drug with a new indication and a new dosage for the treatment of newly-diagnosed chronic phase chronic myeloid leukemia.